PMDA approval: Pentasa Suppositories 1 g
Applicant: Kyorin Pharmaceutical Co., Ltd. Approval date: 2013-03-25. Type: New approval. Active ingredient(s): Mesalazine.
Notes: A drug in a new dosage form indicated for the treatment of ulcerative colitis (excluding severe cases).